PMDA approval: Grtpa Inj. 6,000,000 Grtpa Inj. 12,000,000 Grtpa Inj. 24,000,000 (Mitsubishi Tanabe Pharma Corporation) Activacin for Injection 6,000,000 Activacin for Injection 12,000,000 Activacin for Injection 24,000,000
Applicant: Kyowa Hakko Kirin Co., Ltd. Approval date: 2013-02-28. Type: Partial change. Active ingredient(s): Alteplase (genetical recombination).
Notes: Drugs with a new indication and a new dosage for the improvement of functional impairment in association with acute phase of ischemic cerebrovascular disorder (within 4.5 hours after onset). [Public knowledge-based application after PAFSC's preliminary assessment]